PMDA approval: Voluven Solution for Infusion 6%
Applicant: Fresenius Kabi Japan K.K. Approval date: 2013-03-25. Type: New approval. Active ingredient(s): Hydroxyethylated starch 130000.
Notes: A drug with a new active ingredient indicated for maintenance of circulating blood volume.